PMDA approval: Firazyr Subcutaneous Injection 30 mg Syringe
Applicant: Takeda Pharmaceutical Company Limited. Approval date: 2022-08-24. Type: Partial change. Active ingredient(s): Icatibant acetate.
Notes: A drug with a new additional pediatric dosage indicated for the treatment of acute attacks of hereditary angioedema. [Orphan drug]